PMDA approval: Valixa Tablets 450 mg
Applicant: Mitsubishi Tanabe Pharma Corporation. Approval date: 2009-05-20. Type: Partial change. Active ingredient(s): Valganciclovir hydrochloride.
Notes: A drug with a new additional indication for the treatment of cytomegalovirus infection in organ transplantation (including hematogenic stem cell transplantation), malignant tumor, etc.